PMDA approval: Veltassa 8.4 g Powder for Suspension (Single-dose package)
Applicant: Zeria Pharmaceutical Co., Ltd. Approval date: 2024-09-24. Type: New approval. Active ingredient(s): Patiromer sorbitex calcium.
Notes: A drug with a new active ingredient indicated for the treatment of hyperkalaemia.